PMDA approval: Bavencio Injection 200 mg
Applicant: Merck Biopharma Co., Ltd. Approval date: 2019-12-20. Type: Partial change. Active ingredient(s): Avelumab (genetical recombination).
Notes: A drug with a new indication and a new dosage for the treatment of unresectable or metastatic renal cell carcinoma.